PMDA approval: Cialis Tablets 5 mg Cialis Tablets 10 mg Cialis Tablets 20 mg
Applicant: Eli Lilly Japan K.K. Approval date: 2007-07-31. Type: New approval. Active ingredient(s): Tadalafil.
Notes: Drugs containing a new active ingredient indicated for the treatment of erectile dysfunction (patients unable to obtain or maintain erections sufficient for satisfactory sexual activity).